PMDA approval: Recalbon Tablets 50 mg (Ono Pharmaceutical Co., Ltd.) Bonoteo Tablets 50 mg
Applicant: Astellas Pharma Inc. Approval date: 2011-07-01. Type: New approval. Active ingredient(s): Minodronic acid hydrate.
Notes: Drugs with a new dosage and an additional dosage form indicated for the treatment of osteoporosis.